PMDA approval: Azilva Granules 1% Azilva Tablets 10 mg Azilva Tablets 20 mg Azilva Tablets 40 mg
Applicant: Takeda Pharmaceutical Company Limited. Approval date: 2021-09-27. Type: New approval. Active ingredient(s): Azilsartan.
Notes: Drugs with a new additional pediatric dosage and a drug in an additional dosage form indicated for the treatment of hypertension.